PMDA approval: Humira 40 mg for S.C. Injection Syringe 0.8 mL
Applicant: AbbVie GK. Approval date: 2016-06-20. Type: Partial change. Active ingredient(s): Adalimumab (genetical recombination).
Notes: A drug with a new dosage indicated for the treatment of Crohn's disease.